PMDA approval: Targretin Capsules 75 mg
Applicant: Minophagen Pharmaceutical Co., Ltd. Approval date: 2016-01-22. Type: New approval. Active ingredient(s): Bexarotene.
Notes: A drug with a new active ingredient indicated for the treatment of cutaneous T-cell lymphoma. [Orphan drug]